PMDA approval: Prevymis Tablets 240 mg Prevymis Intravenous Infusion 240 mg Prevymis Granules 20 mg Prevymis Granules 120 mg
Applicant: MSD K.K. Approval date: 2025-03-27. Type: Partial change. Active ingredient(s): Letermovir.
Notes: (1) Drugs with a new pediatric dosage in an additional dosage form indicated for the prophylaxis of cytomegalovirus disease in organ transplantation. (2) Drugs with a new pediatric dosage in an additional dosage form indicated for the prophylaxis of cytomegalovirus disease in allogeneic hematopoietic stem cell transplantation. [Orphan Drug (only for [2])]